PMDA approval: Eslax Intravenous 1%
Applicant: Organon Japan. Approval date: 2007-07-31. Type: New approval. Active ingredient(s): Rocuronium bromide.
Notes: A drug containing a new active ingredient indicated for inducing muscular relaxation during anesthesia and tracheal intubation.